PMDA approval: Mearubik II Subcutaneous Injection
Applicant: The Research Foundation for Microbial Diseases of Osaka University. Approval date: 2024-09-24. Type: New approval. Active ingredient(s): Live attenuated measles virus (Tanabe strain) and live attenuated rubella virus (Matsuura/J16 strain).
Notes: A new combination drug with a new active ingredient indicated for the prevention of measles and rubella.